PMDA approval: Caduet Combination Tablets 1ban Caduet Combination Tablets 2ban Caduet Combination Tablets 3ban Caduet Combination Tablets 4ban
Applicant: Pfizer Japan Inc. Approval date: 2009-07-07. Type: New approval. Active ingredient(s): Amlodipine besilate/atorvastatin calcium hydrate.
Notes: New combination drugs indicated for the treatment of comorbidity of hypertension or angina pectoris and hypercholesterolemia or familial hypercholesterolemia.